PMDA approval: Invega Tablets 3 mg Invega Tablets 6 mg Invega Tablets 9 mg
Applicant: Janssen Pharmaceutical K.K. Approval date: 2010-10-27. Type: New approval. Active ingredient(s): Paliperidone.
Notes: Drugs with a new active ingredient indicated for the treatment of schizophrenia.